PMDA approval: Zykadia Capsules 150 mg
Applicant: Novartis Pharma K.K. Approval date: 2017-09-22. Type: Partial change. Active ingredient(s): Ceritinib.
Notes: A drug with a revised indication for the treatment of unresectable advanced/relapsed ALK fusion gene-positive non-small-cell lung cancer.